PMDA approval: Andembry S.C. Injection 200 mg Pens
Applicant: CSL Behring K.K. Approval date: 2025-02-20. Type: New approval. Active ingredient(s): Garadacimab (genetical recombination).
Notes: A drug with a new active ingredient indicated for the prophylaxis of acute attacks of hereditary angioedema.